PMDA approval: Nuvaxovid Intramuscular Injection 1 mL
Applicant: Takeda Pharmaceutical Company Limited. Approval date: 2024-09-05. Type: New approval. Active ingredient(s): SARS-CoV-2 rS (Omicron JN.1 variant).
Notes: A drug with a new indication, a new dosage, and other characteristics in an additional dosage form for the prevention of disease caused by SARS-CoV-2 infection (COVID-19).